PMDA approval: Sigmart Injection 2 mg Sigmart Injection 12 mg Sigmart Injection 48 mg
Applicant: Chugai Pharmaceutical Co., Ltd. Approval date: 2007-10-19. Type: Partial change. Active ingredient(s): Nicorandil.
Notes: Drugs with a new indication and dosage for the treatment of acute cardiac failure including the acute exacerbation period of chronic cardiac failure.